PMDA approval: Amerge Tablets 2.5 mg
Applicant: GlaxoSmithKline K.K. Approval date: 2008-01-25. Type: New approval. Active ingredient(s): Naratriptan hydrochloride.
Notes: A drug containing a new active ingredient indicated for the treatment of migraine.